PMDA approval: Picoprep Combination Powder
Applicant: Ferring Pharmaceuticals Co.,Ltd. Approval date: 2016-07-04. Type: New approval. Active ingredient(s): Sodium picosulfate hydrate/ Magnesium oxide/ Anhydrous citric acid.
Notes: A new combination drug indicated for bowel cleansing preparation for colonoscopy or colorectal surgery.